PMDA approval: Treakisym Injection 25 mg
Applicant: SymBio Pharmaceuticals Limited. Approval date: 2016-09-28. Type: New approval. Active ingredient(s): Bendamustine hydrochloride.
Notes: A drug with a new additional indication, dosage, and dosage form for the treatment of chronic lymphocytic leukemia. [Orphan drug]